PMDA approval: Primaquine Tablets 15 mg "Sanofi"
Applicant: Sanofi K.K. Approval date: 2016-03-28. Type: New approval. Active ingredient(s): Primaquine phosphate.
Notes: A drug with a new active ingredient indicated for the treatment of malaria caused by Plasmodium vivax and Plasmodium oval.